PMDA approval: Famvir Tab. 250 mg
Applicant: Asahi Kasei Pharma Corporation. Approval date: 2013-02-21. Type: Partial change. Active ingredient(s): Famciclovir.
Notes: A drug with a new additional indication and a new dosage for the treatment of herpes simplex. Drugs with a new additional indication for the treatment of gastritis induced by Helicbacter pylori infection. (9) Pasetocin Capsules 125 Pasetocin Capsules 250 (10) Pasetocin Tablets 250 (Kyowa Hakko Kirin Co., Ltd.) (11) Sawacillin Capsules 125 Sawacillin Capsules 250 (12) Sawacillin Tablets 250 (Astellas Pharma Inc.) (13) Amolin Capsules 125 Amolin Capsules 250 (14) 10% Amolin Fine Granules (Takeda Pharmaceutical Company Limited) (15) Clarith tab. 200 (Taisho Pharmaceutical Co., Ltd.) (16) Klaricid Tablets 200 mg (Abbott Japan Co., Ltd.) (17) Clarithromycin Tab. 200 mg "Mylan" (Mylan Seiyaku Ltd.) (18) Clarithromycin Tablets 200 mg "Takata" (Takata Seiyaku Co., Ltd.) (19) Flagyl Oral Tablet 250 mg (Shionogi & Co., Ltd.) Change Change Change Change Change Change Change Change Change Change Change Change Change Change (21) Lansoprazole/amoxicillin hydrate/metronidazole (20) Lansap 400 Lansap 800 (21) Lampion Pack (Takeda Pharmaceutical Company Limited) Change Change Change